PMDA approval: Miticure House Dust Mite Sublingual Tablets 3,300 JAU Miticure House Dust Mite Sublingual Tablets 10,000 JAU
Applicant: Torii Pharmaceutical Co., Ltd. Approval date: 2018-02-16. Type: Partial change. Active ingredient(s): Dermatophagoides farinae extract, Dermatophagoides pteronyssinus extract.
Notes: Drugs with a new additional pediatric dosage for children aged under 12 years. These drugs comprise allergen immunotherapy for the treatment of house dust mite antigen-induced allergic rhinitis.